PMDA approval: Hemlibra s.c. 30 mg Hemlibra s.c. 60 mg Hemlibra s.c. 90 mg Hemlibra s.c. 105 mg Hemlibra s.c. 150 mg
Applicant: Chugai Pharmaceutical Co., Ltd. Approval date: 2018-12-21. Type: Partial change. Active ingredient(s): Emicizumab (genetical recombination).
Notes: Drugs with a new dosage indicated for the control of bleeding tendency in patients with congenital blood coagulation factor VIII deficiency with blood coagulation factor VIII inhibitors. [Orphan drug]